PMDA approval: EquMet Combination Tablets LD EquMet Combination Tablets HD
Applicant: Novartis Pharma K.K. Approval date: 2015-09-28. Type: New approval. Active ingredient(s): Vildagliptin/Metformin hydrochloride.
Notes: New combination drugs indicated for the treatment of type 2 diabetes mellitus (only when a concomitant use of vildagliptin with metformin hydrochloride is deemed appropriate).